PMDA approval: Ostabalo Subcutaneous Injection Cart 3 mg
Applicant: Teijin Pharma Limited. Approval date: 2021-03-23. Type: New approval. Active ingredient(s): Abaloparatide acetate.
Notes: A drug with a new active ingredient indicated for the treatment of osteoporosis with a high risk of fracture.